PMDA approval: Lynparza Tablets 100 mg Lynparza Tablets 150 mg
Applicant: AstraZeneca K.K. Approval date: 2024-11-22. Type: Partial change. Active ingredient(s): Olaparib.
Notes: Drugs with a new indication and a new dosage for the maintenance therapy of advanced or recurrent endometrial cancer with proficient mismatch repair (pMMR) in patients who have received chemotherapy including treatment with durvalumab (genetical recombination).